PMDA approval: Adsorbed Pandemic Influenza Vaccine (H5N1) "Hokken"
Applicant: The Kitasato Institute. Approval date: 2007-10-19. Type: New approval. Active ingredient(s): Inactivated influenza virus, H5N1 (whole virion).
Notes: A drug containing a new active ingredient indicated for the prevention of pandemic influenza (H5N1). [Orphan drug]